PMDA approval: Graceptor Capsules 0.5 mg Graceptor Capsules 1 mg Graceptor Capsules 5 mg
Applicant: Astellas Pharma Inc. Approval date: 2008-07-16. Type: New approval. Active ingredient(s): Tacrolimus hydrate.
Notes: Drugs in a new dosage form indicated for suppression of organ rejection in renal, liver, heart, lung, and pancreatic transplantation, as well as for suppression of graft rejection and graft versus host disease (GVHD) in bone marrow transplantation.